PMDA approval: Privigen 10% I.V. Drip Infusion 5 g/50 mL Privigen 10% I.V. Drip Infusion 10 g/100 mL Privigen 10% I.V. Drip Infusion 20 g/200 mL
Applicant: CSL Behring K.K. Approval date: 2020-02-21. Type: Partial change. Active ingredient(s): pH4-treated acidic normal human immunoglobulin.
Notes: Drugs with new indications and a new dosage for the treatment of agammaglobulinemia or hypogammaglobulinemia.